PMDA approval: Dopram Injectable 400 mg
Applicant: Kissei Pharmaceutical Co., Ltd. Approval date: 2015-03-20. Type: Partial change. Active ingredient(s): Doxapram hydrochloride hydrate.
Notes: A drug with a new additional indication and a new dosage for the treatment of primary apnoea (apnea of prematurity) in immature or low birth weight infants who have not responded sufficiently to conventional therapies.